PMDA approval: Imovax Polio Subcutaneous Injection
Applicant: Sanofi Pasteur. Approval date: 2012-04-27. Type: New approval. Active ingredient(s): Inactivated poliomyelitis vaccine (Salk vaccine).
Notes: A drug with a new active ingredient indicated for the prevention of acute poliomyelitis. [Expedited review]